PMDA approval: Keytruda Injection 100 mg
Applicant: MSD K.K. Approval date: 2024-05-17. Type: Partial change. Active ingredient(s): Pembrolizumab (genetical recombination).
Notes: A drug with a new indication and a new dosage for the treatment of unresectable biliary tract cancer.